PMDA approval: Lamictal Tablets 2 mg for Children Lamictal Tablets 5 mg for Children Lamictal Tablets 25 mg Lamictal Tablets 100 mg
Applicant: GlaxoSmithKline K.K. Approval date: 2008-10-16. Type: New approval. Active ingredient(s): Lamotrigine.
Notes: Drugs containing a new active ingredient indicated for use as adjunctive treatment with other antiepileptic drugs for partial seizures (including secondary generalized seizures), tonic-clonic seizures, and generalized seizures associated with Lennox-Gastaut syndrome in patients with epilepsy for whom other antiepileptic drugs are not sufficiently effective.